PMDA approval: Lynparza Tablets 100 mg Lynparza Tablets 150 mg
Applicant: AstraZeneca K.K. Approval date: 2023-08-23. Type: Partial change. Active ingredient(s): Olaparib.
Notes: Drugs with a new dosage indicated for the treatment of BRCA mutation-positive, metastatic castration-resistant prostate cancer.